PMDA approval: Adriacin Injection 10 Adriacin Injection 50
Applicant: Kyowa Hakko Kirin Co., Ltd. Approval date: 2014-12-18. Type: Partial change. Active ingredient(s): Doxorubicin hydrochloride.
Notes: Drugs with a revised indication and a new dosage for the relief of symptoms of malignant lymphoma. [Expedited review]